PMDA approval: Tafinlar Capsules 50 mg Tafinlar Capsules 75 mg
Applicant: Novartis Pharma K.K. Approval date: 2018-03-23. Type: Partial change. Active ingredient(s): Dabrafenib mesilate.
Notes: Drugs with a new additional indication and a new dosage indicated for the treatment of unresectable advanced or recurrent BRAF mutation-positive non-small-cell lung cancer. [Orphan drug]